PMDA approval: Gentacin Injection 10 Gentacin Injection 40 Gentacin Injection 60
Applicant: MSD K.K. Approval date: 2013-09-13. Type: Partial change. Active ingredient(s): Gentamicin sulfate.
Notes: Drugs with a new dosage indicated for the treatment of sepsis, secondary infection of trauma, burn, and surgical wound, pneumonia, cystitis, pyelonephritis, peritonitis, otitis media caused by following applicable microorganisms: Gentamicin-sensitive Staphylococcus, Escherichia coli, Klebsiella, Enterobacter, Serratia, Proteus, Morganella morganii, Providencia, Pseudomonas aeruginosa